PMDA approval: Cosopt Ophthalmic Solution
Applicant: Banyu Pharmaceutical Co., Ltd. Approval date: 2010-04-16. Type: New approval. Active ingredient(s): Dorzolamide hydrochloride/timolol maleate.
Notes: A new combination drug indicated for the treatment of glaucoma and ocular hypertension in patients who have not responded sufficiently to other glaucoma drugs.